PMDA approval: Memary Tablets 5 mg Memary Tablets 10 mg Memary Tablets 20 mg
Applicant: Daiichi Sankyo Company, Limited. Approval date: 2011-01-21. Type: New approval. Active ingredient(s): Memantine hydrochloride.
Notes: Drugs with a new active ingredient indicated for inhibition of progression of symptoms of dementia in moderate and severe Alzheimer's dementia.